PMDA approval: Tecentriq Intravenous Infusion 840 mg
Applicant: Chugai Pharmaceutical Co., Ltd. Approval date: 2019-09-20. Type: New approval. Active ingredient(s): Atezolizumab (genetical recombination).
Notes: A drug with a new indication and a new dosage in an additional dosage form indicated for the treatment of PD-L1-positive, hormone receptor-negative and HER2-negative inoperable or recurrent breast cancer.